PMDA approval: (1) Nobelzin Granules 5% (2) Nobelzin Tablets 25 mg (3) Nobelzin Tablets 50 mg
Applicant: Nobelpharma Co., Ltd. Approval date: 2021-01-22. Type: New approval. Active ingredient(s): Zinc acetate hydrate.
Notes: Drugs with a new dosage and a drug in an additional dosage form for the treatment of hypozincemia.